PMDA approval: Prezista Tablets 300 mg
Applicant: Janssen Pharmaceutical K.K. Approval date: 2007-11-22. Type: New approval. Active ingredient(s): Darunavir ethanolate.
Notes: A drug containing a new active ingredient indicated for the treatment of HIV infection. [Orphan drug]